PMDA approval: Fycompa Fine Granules 1% Fycompa Tablets 2 mg Fycompa Tablets 4 mg
Applicant: Eisai Co., Ltd. Approval date: 2020-01-23. Type: New approval. Active ingredient(s): Perampanel hydrate.
Notes: A drug in an additional dosage form and drugs with a revised indication and a new additional pediatric dosage for the treatment of partial seizures (including secondary generalized seizures) in patients with epilepsy.